PMDA approval: Zebiax Lotion 2%
Applicant: Maruho Co., Ltd. Approval date: 2015-09-28. Type: New approval. Active ingredient(s): Ozenoxacin.
Notes: A drug with a new active ingredient indicated for the treatment of superficial skin infections and acne (accompanied by purulent inflammation).